PMDA approval: Pemazyre Tablets 4.5 mg
Applicant: Incyte Biosciences Japan GK. Approval date: 2021-03-23. Type: New approval. Active ingredient(s): Pemigatinib.
Notes: A drug with a new active ingredient indicated for the treatment of unresectable FGFR2 fusion gene-positive biliary tract cancer that has progressed after cancer chemotherapy. [Orphan drug]